PMDA approval: Tracleer pediatric dispersible tablets 32 mg
Applicant: Actelion Pharmaceuticals Japan Ltd. Approval date: 2015-09-28. Type: New approval. Active ingredient(s): Bosentan hydrate.
Notes: A drug with a new indication and a new dosage in a new additional dosage form indicated for the treatment of pulmonary arterial hypertension. [Orphan drug]